PMDA approval: Insulin Aspart BS Injection SoloStar NR "Sanofi" Insulin Aspart BS Injection Cart NR "Sanofi" Insulin Aspart BS Injection 100 I.U./mL NR "Sanofi"
Applicant: Sanofi K.K. Approval date: 2021-03-23. Type: New approval. Active ingredient(s): Insulin aspart (genetical recombination) [insulin aspart biosimilar 1].
Notes: Follow-on biologics indicated for the treatment of diabetes mellitus in cases where insulin therapy is indicated.